PMDA approval: Ibrance Capsules 25 mg Ibrance Capsules 125 mg
Applicant: Pfizer Japan Inc. Approval date: 2017-09-27. Type: New approval. Active ingredient(s): Palbociclib.
Notes: Drugs with a new active ingredient indicated for the treatment of unresectable or recurrent breast cancer.